PMDA approval: Vaxem Hib Suspension Liquid for Injection
Applicant: Takeda Pharmaceutical Company Limited. Approval date: 2016-12-19. Type: Partial change. Active ingredient(s): Haemophilus influenzae type b vaccine adsorbed (mutated diphtheria toxin CRM197 conjugate).
Notes: A drug with a new dosage indicated for the prophylaxis of Haemophilus influenzae type b infections.